PMDA approval: Daichirona for Intramuscular Injection
Applicant: Daiichi Sankyo Company, Limited. Approval date: 2023-08-02. Type: New approval. Active ingredient(s): Ufrenmeran.
Notes: A drug with a new active ingredient indicated for the prevention of disease caused by SARS-CoV-2 infection (COVID-19). [Priority review]